PMDA approval: Farydak Capsules 10 mg Farydak Capsules 15 mg
Applicant: Novartis Pharma K.K. Approval date: 2015-07-03. Type: New approval. Active ingredient(s): Panobinostat lactate.
Notes: Drugs with a new active ingredient indicated for the treatment of relapsed or refractory multiple myeloma. [Orphan drug]